PMDA approval: Thymoglobuline for Intravenous Infusion 25 mg
Applicant: Genzyme Japan K.K. Approval date: 2011-04-22. Type: Partial change. Active ingredient(s): Anti-human thymocyte immunoglobulin, rabbit.
Notes: A drug with a new additional indication and a new dosage for the treatment of acute rejection after renal transplantation. [Expedited review]